PMDA approval: Respia Injection or oral solution 60 mg
Applicant: Nobelpharma Co., Ltd. Approval date: 2014-03-24. Type: New approval. Active ingredient(s): Anhydrous caffeine.
Notes: A drug with a new route of administration indicated for the treatment of primary apnea (apnea of prematurity) in immature or low birth weight infants. [Orphan drug]